PMDA approval: Xtandi Tablets 40 mg Xtandi Tablets 80 mg
Applicant: Astellas Pharma Inc. Approval date: 2020-05-29. Type: Partial change. Active ingredient(s): Enzalutamide.
Notes: Drugs with a new indication for the treatment of metastatic prostate cancer.